PMDA approval: Desalex Tablets 5 mg
Applicant: MSD K.K. Approval date: 2016-09-28. Type: New approval. Active ingredient(s): Desloratadine.
Notes: A drug with a new active ingredient indicated for the treatment of allergic rhinitis, urticaria, and itching associated with skin diseases (eczema/dermatitis, cutaneous pruritus).